PMDA approval: Sprycel Tablets 20 mg Sprycel Tablets 50 mg
Applicant: Bristol-Myers K.K. Approval date: 2009-01-21. Type: New approval. Active ingredient(s): Dasatinib hydrate.
Notes: Drugs containing a new active ingredient indicated for the treatment of 1) imatinib-resistant chronic myelogenous leukemia and 2) recurrent or refractory Philadelphia chromosome-positive acute lymphoblastic leukemia. [Orphan drug]